PMDA approval: Inderal Tablets 10 mg Inderal Tablets 20 mg
Applicant: AstraZeneca K.K. Approval date: 2013-02-28. Type: Partial change. Active ingredient(s): Propranolol hydrochloride.
Notes: Drugs with a new additional indication and a new dosage for the prevention of migraine attack. [Public knowledge-based application after PAFSC's preliminary assessment]